PMDA approval: Hizentra 20% S.C. Injection 1 g/5 mL Hizentra 20% S.C. Injection 2 g/10 mL Hizentra 20% S.C. Injection 4 g/20 mL
Applicant: CSL Behring K.K. Approval date: 2023-06-26. Type: Partial change. Active ingredient(s): Human normal immunoglobulin G (pH4-treated acidic normal human immunoglobulin [subcutaneous injection]).
Notes: Drugs with a new dosage indicated for the treatment of agammaglobulinemia or hypogammaglobulinemia.